PMDA approval: Otezla Tablets 10 mg Otezla Tablets 20 mg Otezla Tablets 30 mg
Applicant: Celgene K.K. Approval date: 2016-12-19. Type: New approval. Active ingredient(s): Apremilast.
Notes: Drugs with a new active ingredient indicated for the treatment of plaque psoriasis in patients who have not responded sufficiently to topical therapy or psoriatic arthritis.